PMDA approval: Azilva Tablets 20 mg Azilva Tablets 40 mg
Applicant: Takeda Pharmaceutical Company Limited. Approval date: 2012-01-18. Type: New approval. Active ingredient(s): Azilsartan.
Notes: Drugs with a new active ingredient indicated for the treatment of hypertension.